PMDA approval: Doxorubicin Hydrochloride for Injection 10 mg “NK” Doxorubicin Hydrochloride for Injection 50 mg “NK”
Applicant: Nippon Kayaku Co., Ltd. Approval date: 2014-12-18. Type: Partial change. Active ingredient(s): Doxorubicin hydrochloride.
Notes: Drugs with a revised indication and a new dosage for the relief of symptoms of malignant lymphoma. [Expedited review]